PMDA approval: Methylene Blue Intravenous Injection 50 mg "Daiichi Sankyo"
Applicant: Daiichi Sankyo Company, Limited. Approval date: 2014-12-26. Type: New approval. Active ingredient(s): Methylthioninium chloride hydrate.
Notes: A drug with a new active ingredient indicated for the treatment of toxic methemoglobinemia.